PMDA approval: Kolbet Tablets 25 mg (Toyama Chemical Co., Ltd.) Careram Tablets 25 mg
Applicant: Eisai Co., Ltd. Approval date: 2012-06-29. Type: New approval. Active ingredient(s): Iguratimod.
Notes: Drugs with a new active ingredient indicated for the treatment of rheumatoid arthritis.